PMDA approval: Mohrus Tape 20 mg Mohrus Tape L 40 mg
Applicant: Hisamitsu Pharmaceutical Co., Inc. Approval date: 2009-11-06. Type: Partial change. Active ingredient(s): Ketoprofen.
Notes: Drugs with a new additional indication for relief of local pain associated with rheumatoid arthritis.